PMDA approval: Afinitor Tablets 2.5 mg Afinitor Tablets 5 mg Afinitor Dispersible Tablets 2 mg Afinitor Dispersible Tablets 3 mg
Applicant: Novartis Pharma K.K. Approval date: 2019-08-22. Type: Partial change. Active ingredient(s): Everolimus.
Notes: Drugs with a new indication and a new dosage for the treatment of tuberous sclerosis complex. [Orphan drug]